PMDA approval: Precedex Intravenous Solution 200 μg "Pfizer" Precedex Intravenous Solution 200 μg/50 mL Syringe "Pfizer" (Pfizer Japan Inc.) Precedex Intravenous Solution 200 μg "Maruishi" Precedex Intravenous Solution 200 μg/50 mL Syringe "Maruishi"
Applicant: Maruishi Pharmaceutical Co., Ltd. Approval date: 2018-11-29. Type: Partial change. Active ingredient(s): Dexmedetomidine hydrochloride.
Notes: Drugs with a new additional pediatric dosage indicated for sedation during artificial respiration and after weaning in patients in intensive care.